PMDA approval: Tepmetko Tablets 250 mg
Applicant: Merck Biopharma Co., Ltd. Approval date: 2020-03-25. Type: New approval. Active ingredient(s): Tepotinib hydrochloride hydrate.
Notes: A drug with a new active ingredient indicated for the treatment of unresectable advanced or recurrent MET exon 14 (METex14) skipping mutation-positive non-small cell lung cancer. [SAKIGAKE designation] [Orphan drug]